PMDA approval: Ciclosporin Cap. 10 mg "Mylan" Ciclosporin Cap. 25 mg "Mylan" Ciclosporin Cap. 50 mg "Mylan" Ciclosporin Fine Granules 17% "Mylan"
Applicant: Mylan Seiyaku Ltd. Approval date: 2013-03-25. Type: Partial change. Active ingredient(s): Ciclosporin.
Notes: Drugs with a new indication and a new dosage for the treatment of non-Behcet's, non-infectious uveitis (active non-infectious uveitis in the intermediate or posterior area in patients who have not responded sufficiently to conventional treatments and may involve deterioration of visual acuity). [Public knowledge-based application after PAFSC's preliminary assessment]